PMDA approval: Eybelis Ophthalmic Solution 0.002%
Applicant: Santen Pharmaceutical Co., Ltd. Approval date: 2018-09-21. Type: New approval. Active ingredient(s): Omidenepag isopropyl.
Notes: A drug with a new active ingredient indicated for the treatment of glaucoma and ocular hypertension.